PMDA approval: Xospata Tablets 40 mg
Applicant: Astellas Pharma Inc. Approval date: 2018-09-21. Type: New approval. Active ingredient(s): Gilteritinib fumarate.
Notes: A drug with a new active ingredient indicated for the treatment of relapsed or refractory FLT3 mutation-positive acute myeloid leukemia. [Orphan drug, SAKIGAKE designation]